PMDA approval: Rituximab BS Intravenous Infusion 100 mg [KHK] Rituximab BS Intravenous Infusion 500 mg [KHK]
Applicant: Sandoz K.K. Approval date: 2017-09-27. Type: New approval. Active ingredient(s): Rituximab (genetical recombination) [Rituximab biosimilar 1].
Notes: Follow-on biologics indicated for the treatment of CD20-positive B-cell non-Hodgkin's lymphoma, CD20-positive B-cell lymphoproliferative disorder associated with immunosuppression, Wegener's granulomatosis, and microscopic polyangiitis.